PMDA approval: Ezharmia Tablets 50 mg Ezharmia Tablets 100 mg
Applicant: Daiichi Sankyo Company, Limited. Approval date: 2022-09-26. Type: New approval. Active ingredient(s): Valemetostat tosilate.
Notes: Drugs with a new active ingredient indicated for the treatment of relapsed or refractory adult T-cell leukemia/lymphoma. [Orphan drug]